PMDA approval: Fibrogammin P I.V. Injection
Applicant: CSL Behring K.K. Approval date: 2013-09-13. Type: Partial change. Active ingredient(s): Lyophilized human blood coagulation factor XIII concentrate.
Notes: A drug with a new additional indication for the treatment of bleeding tendency caused by acquired blood coagulation factor XIII deficiency. [Public knowledge-based application after PAFSC's preliminary assessment]